PMDA approval: Midafresa Injection 0.1%
Applicant: Alfresa Pharma Corporation. Approval date: 2021-09-27. Type: Partial change. Active ingredient(s): Midazolam.
Notes: A drug with a new dosage indicated for the treatment of status epilepticus.